PMDA approval: Livtencity Tablets 200 mg
Applicant: Takeda Pharmaceutical Company Limited. Approval date: 2024-06-24. Type: New approval. Active ingredient(s): Maribavir.
Notes: A drug with a new active ingredient indicated for the treatment of cytomegalovirus disease in organ transplantation (including hematopoietic stem cell transplantation) that is refractory to conventional anti-cytomegalovirus therapy. [Orphan drug]